PMDA approval: Diovan Tablets 20 mg Diovan Tablets 40 mg Diovan Tablets 80 mg Diovan Tablets 160 mg
Applicant: Novartis Pharma K.K. Approval date: 2012-08-24. Type: Partial change. Active ingredient(s): Valsartan.
Notes: Drugs with a new additional pediatric dosage. These drugs are indicated for the treatment of hypertension. [Public knowledge-based application after PAFSC's preliminary assessment]